PMDA approval: Duodopa Enteral Combination Solution
Applicant: AbbVie GK. Approval date: 2016-07-04. Type: New approval. Active ingredient(s): Levodopa/ Carbidopa hydrate.
Notes: A new combination drug indicated for the improvement of wearing-off phenomenon in the symptoms of Parkinson's disease in patients who have not responded sufficiently to conventional levodopa-containing drug therapies. [Orphan drug]